PMDA approval: Entyvio for I.V. Infusion 300 mg
Applicant: Takeda Pharmaceutical Company Limited. Approval date: 2018-07-02. Type: New approval. Active ingredient(s): Vedolizumab (genetical recombination).
Notes: A drug with a new active ingredient indicated for the treatment and maintenance therapy of moderate to severe ulcerative colitis (for use only in patients who have not sufficiently responded to conventional treatments).